PMDA approval: Biopten Granules 2.5%
Applicant: Asubio Pharma Co., Ltd. Approval date: 2008-07-16. Type: Partial change. Active ingredient(s): Sapropterin hydrochloride.
Notes: A drug with a new indication and a new dosage for reduction of serum phenylalanine levels in patients with hyperphenylalaninemia (tetrahydrobiopterin-responsive hyperphenylalaninemia) caused by tetrahydrobiopterin-responsive phenylalanine hydroxylase deficiency. [Orphan drug]